PMDA approval: Kenketsu Venoglobulin IH 5% I.V. 2.5 g/50 mL
Applicant: Benesis Corporation. Approval date: 2010-10-27. Type: Partial change. Active ingredient(s): Polyethylene glycol treated human normal immunoglobulin.
Notes: A drug with a new additional indication and a new dosage for the improvement of muscular weakness in polymyositis/dermatomyositis (for use only when steroids are not sufficiently effective). [Orphan drug]